PMDA approval: Tecentriq Intravenous Infusion 840 mg Tecentriq Intravenous Infusion 1200 mg
Applicant: Chugai Pharmaceutical Co., Ltd. Approval date: 2025-09-19. Type: Partial change. Active ingredient(s): Atezolizumab (genetical recombination).
Notes: Drugs with a new dosage indicated for the treatment of following diseases: ・Unresectable advanced or recurrent non-small cell lung cancer ・Postoperative adjuvant treatment for PD-L1 positive non-small cell lung cancer ・PD-L1 positive, hormone receptor (HR)-negative, and human epidermal growth factor receptor 2 (HER2)-negative inoperable or recurrent breast cancer ・Extensive-stage small-cell lung cancer